PMDA approval: Erbitux Injection 100 mg
Applicant: Merck Biopharma Co., Ltd. Approval date: 2022-09-26. Type: Partial change. Active ingredient(s): Cetuximab (genetical recombination).
Notes: A drug with a new additional dosage indicated for the treatment of unresectable, advanced or recurrent colon or rectal cancer with wild-type RAS and head and neck cancer. [Public knowledge-based application]